PMDA approval: Epkinly Subcutaneous Injection 4 mg Epkinly Subcutaneous Injection 48 mg
Applicant: Genmab K.K. Approval date: 2025-02-20. Type: Partial change. Active ingredient(s): Epcoritamab (genetical recombination).
Notes: Drugs with a new dosage indicated for the treatment of relapsed or refractory follicular lymphoma (Grade 1 to Grade 3 A).